PMDA approval: Fastic Tablets 30 Fastic Tablets 90 (Ajinomoto Co., Inc.) Starsis Tablets 30 mg Starsis Tablets 90 mg
Applicant: Astellas Pharma Inc. Approval date: 2007-11-13. Type: Partial change. Active ingredient(s): Nateglinide.
Notes: Drugs with a new indication for reducing glycemic excursions in patients with type 2 diabetes mellitus (in the case where therapy with biguanides in addition to diet and exercise therapy is not sufficiently effective).